PMDA approval: Nubeqa Tablets 300 mg
Applicant: Bayer Yakuhin, Ltd. Approval date: 2020-01-23. Type: New approval. Active ingredient(s): Darolutamide.
Notes: A drug with a new active ingredient indicated for the treatment of non-metastatic, castration-resistant prostate cancer.